PMDA approval: Zagallo Capsules 0.1 mg Zagallo Capsules 0.5 mg
Applicant: GlaxoSmithKline K.K. Approval date: 2015-09-28. Type: New approval. Active ingredient(s): Dutasteride.
Notes: A drug with a new indication and a new dosage in an additional dosage form, and a drug with a new indication and a new dosage, indicated for the treatment of the male pattern hair loss (androgenetic alopecia) in men.